PMDA approval: Humalog Cart Humalog Miriopen
Applicant: Eli Lilly Japan K.K. Approval date: 2016-12-19. Type: Partial change. Active ingredient(s): Insulin lispro (genetical recombination).
Notes: Drugs with a new dosage indicated for the treatment of diabetes mellitus in cases where insulin therapy is indicated.